PMDA approval: Entyvio Pens for S.C. Injection 108 mg Entyvio Syringes for S.C. Injection 108 mg
Applicant: Takeda Pharmaceutical Company Limited. Approval date: 2023-03-27. Type: New approval. Active ingredient(s): Vedolizumab (genetical recombination).
Notes: Drugs with a new route of administration indicated for the maintenance therapy of moderate to severe ulcerative colitis (for use only in patients who have not sufficiently responded to conventional treatments).